PMDA approval: Asacol Tablets 400 mg
Applicant: Zeria Pharmaceutical Co., Ltd. Approval date: 2009-10-16. Type: New approval. Active ingredient(s): Mesalazine.
Notes: A drug in a new dosage form and with a new dosage indicated for the treatment of ulcerative colitis (excluding severe case).